PMDA approval: Feburic Tablets 10 mg Feburic Tablets 20 mg Feburic Tablets 40 mg
Applicant: Teijin Pharma Limited. Approval date: 2016-05-23. Type: Partial change. Active ingredient(s): Febuxostat.
Notes: Drugs with a new additional indication and a new dosage for the treatment of hyperuricemia in patients receiving cancer chemotherapy.